PMDA approval: Nuvaxovid Intramuscular Injection
Applicant: Takeda Pharmaceutical Company Limited. Approval date: 2022-04-19. Type: New approval. Active ingredient(s): Recombinant coronavirus (SARS-CoV-2) vaccine.
Notes: A drug with a new active ingredient indicated for the prevention of disease caused by SARS-CoV-2 infection (COVID-19). [Priority review]